PMDA approval: Enalart Fine Gran. 1% Enalart Tab. 2.5 mg Enalart Tab. 5 mg Enalart Tab. 10 mg
Applicant: Kyowa Pharmaceutical Industry Co., Ltd. Approval date: 2012-06-22. Type: Partial change. Active ingredient(s): Enalapril maleate.
Notes: Drugs with a new additional pediatric dosage. These drugs are indicated for the treatment of hypertension. [Public knowledge-based application after PAFSC's preliminary assessment]